PMDA approval: Remicade for I.V. Infusion 100
Applicant: Mitsubishi Tanabe Pharma Corporation. Approval date: 2015-12-21. Type: Partial change. Active ingredient(s): Infliximab (genetical recombination).
Notes: A drug with a new additional indication and a new dosage for the treatment of acute-phase Kawasaki's disease in patients who have not responded sufficiently to conventional therapies. [Orphan drug]